PMDA approval: Steboronine 9000 mg/300 mL for infusion
Applicant: Stella Pharma Corporation. Approval date: 2020-03-25. Type: New approval. Active ingredient(s): Borofalan (10B).
Notes: A drug with a new active ingredient indicated for the treatment of locally unresectable recurrent or unresectable advanced head and neck cancer. [SAKIGAKE designation]